PMDA approval: Ustekinumab BS 45 mg Syringe for S.C. Injection “F”
Applicant: Fuji Pharma Co., Ltd. Approval date: 2023-09-25. Type: New approval. Active ingredient(s): Ustekinumab (genetical recombination) [ustekinumab biosimilar 1].
Notes: A biosimilar indicated for the treatment of the following disease in patients who have not sufficiently responded to conventional therapies: plaque psoriasis and psoriatic arthritis